PMDA approval: Sarclisa 100 mg I.V. Infusion Sarclisa 500 mg I.V. Infusion
Applicant: Sanofi K.K. Approval date: 2020-06-29. Type: New approval. Active ingredient(s): Isatuximab (genetical recombination).
Notes: Drugs with a new active ingredient indicated for the treatment of relapsed or refractory multiple myeloma.